PMDA approval: Inlyta Tablets 1 mg Inlyta Tablets 5 mg
Applicant: Pfizer Japan Inc. Approval date: 2012-06-29. Type: New approval. Active ingredient(s): Axitinib.
Notes: Drugs with a new active ingredient indicated for the treatment of unresectable or metastatic renal cell carcinoma.